PMDA approval: Prevenar 13 Suspension Liquid for Injection
Applicant: Pfizer Japan Inc. Approval date: 2020-05-29. Type: Partial change. Active ingredient(s): Pneumococcal 13-valent conjugate vaccine adsorbed (mutated diphtheria CRM197 conjugate).
Notes: A drug with a new indication for the prophylaxis of pneumococcal disease (serotypes 1, 3, 4, 5, 6A, 6B, 7F, 9V, 14, 18C, 19A, 19F and 23F) in individuals who are considered to be at high risk of pneumococcal disease.